PMDA approval: Meiact MS Fine Granules 10% for Pediatric
Applicant: Meiji Seika Pharma Co., Ltd. Approval date: 2012-06-22. Type: Partial change. Active ingredient(s): Cefditoren pivoxil.
Notes: A drug with a new additional dosage for the treatment of pneumonia, otitis media, and sinusitis.